PMDA approval: Melatobel Granules 0.2% for Pediatric
Applicant: Nobelpharma Co., Ltd. Approval date: 2020-03-25. Type: New approval. Active ingredient(s): Melatonin.
Notes: A drug with a new active ingredient indicated for the improvement of difficulty falling asleep associated with pediatric neurodevelopmental disorder.